PMDA approval: Exal for Inj. 10 mg
Applicant: Nippon Kayaku Co., Ltd. Approval date: 2011-03-10. Type: Partial change. Active ingredient(s): Vinblastine sulfate.
Notes: A drug with a new additional indication and a new dosage for the treatment of Langerhans cell histiocytosis. [Public knowledge-based application after preliminary assessment by the Pharmaceutical Affairs and Food Sanitation Council (PAFSC)]